PMDA approval: Opdivo Intravenous Infusion 20 mg Opdivo Intravenous Infusion 100 mg
Applicant: Ono Pharmaceutical Co., Ltd. Approval date: 2016-08-26. Type: Partial change. Active ingredient(s): Nivolumab (genetical recombination).
Notes: Drugs with a new additional indication for the treatment of unresectable or metastatic renal cell carcinoma. [Priority review]